PMDA approval: Elplat for Injection 100 mg Elplat I.V. Infusion Solution 50 mg Elplat I.V. Infusion Solution 100 mg
Applicant: Yakult Honsha Co., Ltd. Approval date: 2009-09-18. Type: Partial change. Active ingredient(s): Oxaliplatin.
Notes: Drugs with a new additional indication and a new additional dosage and administration for combination therapy with other anticancer drugs (XELOX + BV regimen) for advanced or recurrent colorectal cancer not suited for curative resection. 87 Elplat for Injection 50 mg (Yakult Honsha Co., Ltd.) Change